PMDA approval: Mepsevii Intravenous Infusion 10 mg
Applicant: Amicus Therapeutics, Inc. Approval date: 2022-01-20. Type: New approval. Active ingredient(s): Vestronidase alfa (genetical recombination).
Notes: A drug with a new active ingredient indicated for the treatment of mucopolysaccharidosis type VII. [Orphan drug]